PMDA approval: Victoza Subcutaneous Injection 18 mg
Applicant: Novo Nordisk Pharma Ltd. Approval date: 2014-08-29. Type: Partial change. Active ingredient(s): Liraglutide (genetical recombination).
Notes: A drug with a new indication for the treatment of type 2 diabetes mellitus.